PMDA approval: Clozaril Tablets 25 mg Clozaril Tablets 100 mg
Applicant: Novartis Pharma K.K. Approval date: 2009-04-22. Type: New approval. Active ingredient(s): Clozapine.
Notes: Drugs with a new active ingredient indicated for the treatment of treatment-resistant schizophrenia.